PMDA approval: Gammagard for Intravenous Injection 2.5 g
Applicant: Baxter Limited. Approval date: 2010-05-13. Type: Partial change. Active ingredient(s): Freeze-dried ion-exchange-resin treated human normal immunoglobulin.
Notes: A drug with a new dosage indicated for the treatment of hypogammaglobulinemia or agammaglobulinemia.